PMDA approval: Bonalon Bag for I.V. Infusion 900 μg
Applicant: Teijin Pharma Limited. Approval date: 2012-01-18. Type: New approval. Active ingredient(s): Alendronate sodium hydrate.
Notes: A drug with a new dosage in a new additional dosage form indicated for the treatment of osteoporosis (not in the reexamination period).